PMDA approval: Meropen for Intravenous Drip Infusion Vial 0.25 g Meropen for Intravenous Drip Infusion Vial 0.5 g Meropen for Intravenous Drip Infusion Kit 0.5 g
Applicant: Dainippon Sumitomo Pharma Co., Ltd. Approval date: 2013-12-20. Type: Partial change. Active ingredient(s): Meropenem hydrate.
Notes: Drugs with a new dosage (the daily dose has been changed) in patients with purulent meningitis.